PMDA approval: Tetrabik Subcutaneous Injection Syringe
Applicant: The Research Foundation for Microbial Diseases of Osaka University. Approval date: 2012-07-27. Type: New approval. Active ingredient(s): Adsorbed diphtheria-purified pertussis-tetanus-inactivated polio (Sabin strain) combined vaccine.
Notes: A drug with a new active ingredient indicated for the prevention of pertussis, diphtheria, tetanus, and acute poliomyelitis.